PMDA approval: Vancomycin Hydrochloride for Intravenous Drip Infusion 0.5 g
Applicant: Shionogi & Co., Ltd. Approval date: 2014-05-23. Type: Partial change. Active ingredient(s): Vancomycin hydrochloride.
Notes: A drug with new additional indications for the treatment of: (1) sepsis, infective endocarditis, secondary infection of trauma, burn or surgical wounds, osteomyelitis, arthritis, peritonitis and purulent meningitis caused by vancomycin-sensitive methicillin-resistant coagulase-negative Staphylococcus (MRCNS); or (2) febrile neutropenia which is suspected of MRSA or MRCNS infection. [Public knowledge-based application after PAFSC's preliminary assessment]